PMDA approval: Freeze-dried Smallpox Vaccine Prepared in Cell Culture LC16 “KMB”
Applicant: KM Biologics Co., Ltd. Approval date: 2022-08-02. Type: Partial change. Active ingredient(s): Live vaccinia virus (LC16m8 strain).
Notes: A drug with a new indication for the prevention of monkeypox.